PMDA approval: Truqap Tablets 160 mg Truqap Tablets 200 mg
Applicant: AstraZeneca K.K. Approval date: 2024-03-26. Type: New approval. Active ingredient(s): Capivasertib.
Notes: Drugs with a new active ingredient indicated for the treatment of hormone receptor (HR)-positive and human epidermal growth factor receptor 2 (HER2)-negative inoperable or recurrent breast cancer with PIK3CA, AKT1, or PTEN mutation that has progressed after endocrine therapy.